PMDA approval: Silgard9 Aqueous Suspension for Intramuscular Injection Syringes
Applicant: MSD K.K. Approval date: 2020-07-21. Type: New approval. Active ingredient(s): Recombinant adsorbed 9-valent human papillomavirus virus-like particle vaccine (yeast origin).
Notes: A drug with a new active ingredient indicated for the prevention of the following diseases caused by infection with human papillomavirus types 6, 11, 16, 18, 31, 33, 45, 52, and 58: • Cervical cancer (squamous cell carcinoma and adenosquamous carcinoma) and its precursor lesions (cervical intraepithelial neoplasia [CIN] 1, 2, and 3, and adenocarcinoma in situ [AIS]) • Vulvar intraepithelial neoplasia (VIN) 1, 2, and 3, and vaginal intraepithelial noeplasia (VaIN) 1, 2, and 3 • Condyloma acuminatum